PMDA approval: Cetrotide for Injection 0.25 mg
Applicant: Nippon Kayaku Co., Ltd. Approval date: 2022-09-16. Type: Partial change. Active ingredient(s): Cetrorelix acetate.
Notes: A drug with a new dosage indicated for the prevention of premature ovulation during controlled ovarian stimulation. [Public knowledge-based application after PAFSC's preliminary assessment]